PMDA approval: Regtect Tablets 333 mg
Applicant: Nippon Shinyaku Co., Ltd. Approval date: 2013-03-25. Type: New approval. Active ingredient(s): Acamprosate calcium.
Notes: A drug with a new active ingredient indicated for use as an aid for maintenance of abstinence in patients with alcohol dependence.